PMDA approval: Qalsody Intrathecal Injection 100 mg
Applicant: Biogen Japan Ltd. Approval date: 2024-12-27. Type: New approval. Active ingredient(s): Tofersen.
Notes: A drug with a new active ingredient indicated for slowing the progression of functional impairment in patients with amyotrophic lateral sclerosis with SOD1 mutation. [Orphan drug]